PMDA approval: Mitchga Syringes 60 mg
Applicant: Maruho Co., Ltd. Approval date: 2022-03-28. Type: New approval. Active ingredient(s): Nemolizumab (genetical recombination).
Notes: A drug with a new active ingredient indicated for the treatment of pruritus associated with atopic dermatitis (for use only in patients who have not responded sufficiently to conventional treatments).